PMDA approval: Modiodal Tablets 100 mg
Applicant: Alfresa Pharma Corporation. Approval date: 2020-02-21. Type: Partial change. Active ingredient(s): Modafinil.
Notes: A drug with a new indication for the treatment of excessive daytime sleepiness associated with idiopathic hypersomnia. [Orphan drug]